PMDA approval: Radicut Oral Suspension 2.1%
Applicant: Mitsubishi Tanabe Pharma Corporation. Approval date: 2022-12-23. Type: New approval. Active ingredient(s): Edaravone.
Notes: A drug with a new route of administration indicated for delaying the functional disorder in patients with amyotrophic lateral sclerosis (ALS).